PMDA approval: Jivi for iv injection 500 Jivi for iv injection 1000 Jivi for iv injection 2000 Jivi for iv injection 3000
Applicant: Bayer Yakuhin, Ltd. Approval date: 2025-09-19. Type: Partial change. Active ingredient(s): Damoctocog alfa pegol (genetical recombination).
Notes: Drugs with a new additional pediatric dosage indicated for the control of bleeding tendency in patients with blood coagulation factor VIII deficiency.